PMDA approval: Xarelto Tablets 10 mg Xarelto Fine Granules 10 mg Xarelto OD Tablets 10 mg Xarelto Dry Syrup for Pediatric 51.7 mg Xarelto Dry Syrup for Pediatric 103.4 mg Xarelto Tablets 2.5 mg
Applicant: Bayer Yakuhin, Ltd. Approval date: 2023-11-24. Type: Partial change. Active ingredient(s): Rivaroxaban.
Notes: Drugs with a new indication and a new dosage for the inhibition of thrombus/embolization formation in patients after Fontan surgery.